PMDA approval: Benlysta for I.V. Infusion 120 mg Benlysta for I.V. Infusion 400 mg Benlysta for S.C. Injection 200 mg Autoinjector Benlysta for S.C. Injection 200 mg Syringe
Applicant: GlaxoSmithKline K.K. Approval date: 2017-09-27. Type: New approval. Active ingredient(s): Belimumab (genetical recombination).
Notes: Drugs with a new active ingredient indicated for the treatment of systemic lupus erythematosus in patients who have not responded sufficiently to conventional treatments.